PMDA approval: Adynovate Intravenous 250 Adynovate Intravenous 500 Adynovate Intravenous 1000 Adynovate Intravenous 2000
Applicant: Baxter Limited. Approval date: 2016-03-28. Type: New approval. Active ingredient(s): Rurioctocog alfa pegol (genetical recombination).
Notes: Drugs with a new active ingredient indicated for the control of bleeding tendency in patients with blood coagulation factor VIII deficiency.